PMDA approval: Denotas Chewable Combination Tablets
Applicant: Nitto Pharmaceutical Industries, Ltd. Approval date: 2013-02-28. Type: New approval. Active ingredient(s): Precipitated calcium carbonate/cholecalciferol/ magnesium carbonate.
Notes: A combination prescription drug with a similar formulation indicated for the treatment and prevention of hypocalcemia in association with administration of RANKL inhibitors (e.g. denosumab [genetical recombination]) [Expedited review]